PMDA approval: Ninlaro Capsules 2.3 mg Ninlaro Capsules 3 mg Ninlaro Capsules 4 mg
Applicant: Takeda Pharmaceutical Company Limited. Approval date: 2021-05-27. Type: Partial change. Active ingredient(s): Ixazomib citrate.
Notes: Drugs with a new indication for the maintenance therapy of multiple myeloma.